PMDA approval: Voranigo Tablets 10 mg Voranigo Tablets 40 mg
Applicant: Nihon Servier Co., Ltd. Approval date: 2025-09-19. Type: New approval. Active ingredient(s): Vorasidenib citric acid hydrate.
Notes: Drugs with a new active ingredient indicated for the treatment of IDH1 or IDH2 gene mutation-positive glioma. [Orphan drug]